PMDA approval: Dupixent 300 mg Syringe for S.C. Injection Dupixent 300 mg Pen for S.C. Injection
Applicant: Sanofi K.K. Approval date: 2025-03-27. Type: Partial change. Active ingredient(s): Dupilumab (genetical recombination).
Notes: Drugs with a new indication and a new dosage for the treatment of chronic obstructive pulmonary disease (for use only in patients who have not responded sufficiently to conventional treatments).